PMDA approval: (1) Omvoh Intravenous Infusion 300 mg (2) Omvoh Subcutaneous Injection 100 mg Autoinjectors (3) Omvoh Subcutaneous Injection 100 mg Syringes
Applicant: Eli Lilly Japan K.K. Approval date: 2023-03-27. Type: New approval. Active ingredient(s): Mirikizumab (genetical recombination).
Notes: (1) A drug with a new active ingredient indicated for the remission induction therapy of moderate to severe ulcerative colitis (for use only in patients who have not sufficiently responded to conventional treatments). (2) (3) Drugs with a new active ingredient indicated for the maintenance therapy of moderate to severe ulcerative colitis (for use only in patients who have not sufficiently responded to conventional treatments).